PMDA approval: Tapros Ophthalmic Solution 0.0015 ％
Applicant: Santen Pharmaceutical Co., Ltd. Approval date: 2008-10-16. Type: New approval. Active ingredient(s): Tafluprost.
Notes: A drug containing a new active ingredient indicated for the treatment of glaucoma and ocular hypertension.